PMDA approval: Femara Tablets 2.5 mg
Applicant: Novartis Pharma K.K. Approval date: 2022-02-25. Type: Partial change. Active ingredient(s): Letrozole.
Notes: A drug with a new indication and a new dosage for controlled ovarian stimulation in assisted reproductive technology. [Public knowledge-based application]